PMDA approval: Revlimid Capsules 2.5 mg Revlimid Capsules 5 mg
Applicant: Celgene K.K. Approval date: 2015-12-21. Type: Partial change. Active ingredient(s): Lenalidomide hydrate.
Notes: Drugs with a revised indication for the treatment of multiple myeloma.